PMDA approval: Arexvy Intramuscular Injection
Applicant: GlaxoSmithKline K.K. Approval date: 2023-09-25. Type: New approval. Active ingredient(s): Recombinant respiratory syncytial virus PreF3 antigen.
Notes: A drug with a new active ingredient indicated for the prevention of disease caused by RS virus infection.